PMDA approval: Symtuza Combination Tablets
Applicant: Janssen Pharmaceutical K.K. Approval date: 2019-06-18. Type: New approval. Active ingredient(s): Darunavir ethanolate/Cobicistat /Emtricitabine/Tenofovir alafenamide fumarate.
Notes: A new combination drug indicated for the treatment of HIV-1 infection. [Orphan drug]